PMDA approval: Kenketu Glovenin-I for I.V. Injection 2500 mg
Applicant: Nihon Pharmaceutical Co., Ltd. Approval date: 2010-05-13. Type: Partial change. Active ingredient(s): Freeze-dried polyethylene glycol treated human normal immunoglobulin.
Notes: A drug with a new dosage indicated for the treatment of hypogammaglobulinemia or agammaglobulinemia.